PMDA approval: Tapcom Combination Ophthalmic Solution
Applicant: Santen Pharmaceutical Co., Ltd. Approval date: 2013-09-20. Type: New approval. Active ingredient(s): (1) Tafluprost (2) Timolol maleate.
Notes: A new combination drug indicated for the treatment of glaucoma and ocular hypertension.